PMDA approval: Alaglio Granules sachets1.5 g
Applicant: SBI Pharmaceuticals Co., Ltd. Approval date: 2017-09-27. Type: New approval. Active ingredient(s): Aminolevulinic acid hydrochloride.
Notes: A drug with a new additional indication and a new dosage in an additional dosage form for the visualization of tumor tissues of the non-muscle invasive bladder cancer in transurethral resection of bladder tumor. [Orphan drug]